PMDA approval: Randa Inj. 10 mg/20 mL Randa Inj. 25 mg/50 mL Randa Inj. 50 mg/100 mL (Nippon Kayaku Co., Ltd.) Briplatin Injection 10 mg Briplatin Injection 25 mg Briplatin Injection 50 mg (Bristol-Myers K.K.) Cisplatin Injection 10 mg 'Nichi-iko' Cisplatin Injection 25 mg 'Nichi-iko' Cisplatin Injection 50 mg 'Nichi-iko' (Nichi-Iko Pharmaceutical Co., Ltd.) Cisplatin for I.V. Infusion 10 mg 'Maruko' Cisplatin for I.V. Infusion 25 mg 'Maruko' Cisplatin for I.V. Infusion 50 mg 'Maruko' (Nichi-Iko Pharmaceutical Co., Ltd.) Platosin Injection 10 Platosin Injection 25 Platosin Injection 50
Applicant: Pfizer Japan Inc. Approval date: 2012-02-22. Type: Partial change. Active ingredient(s): Cisplatin.
Notes: Drugs with a new additional indication and a new dosage for the treatment of biliary tract cancer. [Public knowledge-based application after PAFSC's preliminary assessment]